PMDA approval: Bylvay Granules 200 µg Bylvay Granules 600 µg
Applicant: Ipsen Co., Ltd. Approval date: 2025-09-19. Type: New approval. Active ingredient(s): Odevixibat hydrate.
Notes: Drugs with a new active ingredient indicated for the treatment of pruritus in patients with progressive familial intrahepatic cholestasis. [Orphan drug]